PMDA approval: Adynovate Intravenous 250 Adynovate Intravenous 500 Adynovate Intravenous 1000 Adynovate Intravenous 2000
Applicant: Baxalta Japan Limited. Approval date: 2017-11-30. Type: Partial change. Active ingredient(s): Rurioctocog alfa pegol (genetical recombination).
Notes: Drugs with a new additional pediatric dosage indicated for the control of bleeding tendency in patients with blood coagulation factor VIII deficiency.